PMDA approval: Idelvion I.V. Injection 250 Idelvion I.V. Injection 500 Idelvion I.V. Injection 1000 Idelvion I.V. Injection 2000 Idelvion I.V. Injection 3500
Applicant: CSL Behring K.K. Approval date: 2020-08-21. Type: Partial change. Active ingredient(s): Albutrepenonacog alfa (genetical recombination).
Notes: Drugs with a new dosage indicated for the control of bleeding tendency in patients with blood coagulation factor IX deficiency.